PMDA approval: (1) Simponi Subcutaneous Injection 50 mg Syringe (2) Simponi Subcutaneous Injection 100 mg Syringe
Applicant: Janssen Pharmaceutical K.K. Approval date: 2017-03-30. Type: Partial change. Active ingredient(s): Golimumab (genetical recombination).
Notes: Drugs with a new additional indication and a new dosage, in a new additional dosage form for improvement and maintenance of moderate to severe ulcerative colitis (for use only in patients who have not responded sufficiently to conventional treatments).